PMDA approval: Potassium Iodide Pills 50 mg "Nichi-iko"
Applicant: Nichi-Iko Pharmaceutical Co., Ltd. Approval date: 2013-04-30. Type: Partial change. Active ingredient(s): Potassium iodide.
Notes: A drug with a new additional indication and a new dosage for the prevention and reduction of internal exposure to radioactive iodine in the thyroid gland. [Expedited review]